PMDA approval: Zetia Tablets 10 mg
Applicant: Schering-plough K.K. Approval date: 2007-04-18. Type: New approval. Active ingredient(s): Ezetimibe.
Notes: A drug containing a new active ingredient indicated for the inhibition of cholesterol absorption.